PMDA approval: Ecard Combination Tablets LD Ecard Combination Tablets HD
Applicant: Takeda Pharmaceutical Company Limited. Approval date: 2009-01-21. Type: New approval. Active ingredient(s): Candesartan cilexetil/hydrochlorothiazide.
Notes: New combination drugs indicated for the treatment of hypertension.